PMDA approval: Rethio 100 mg for Intravenous Infusion
Applicant: Sumitomo Dainippon Pharma Co., Ltd. Approval date: 2019-03-26. Type: New approval. Active ingredient(s): Thiotepa.
Notes: A drug with a new active ingredient indicated for the conditioning treatment of solid tumors in pediatric patients prior to autologous hematopoietic stem-cell transplantation. [Expedited review]